PMDA approval: Julina Tablets 0.5 mg
Applicant: Bayer Yakuhin, Ltd. Approval date: 2008-04-16. Type: New approval. Active ingredient(s): Estradiol.
Notes: A drug with a new route of administration indicated for the treatment of vasomotor symptoms (hot flushes and sweating) and vaginal atrophy symptoms associated with climacteric disturbance and ovarian deficiency symptoms.